PMDA approval: Zentacort Capsules 3 mg
Applicant: Zeria Pharmaceutical Co., Ltd. Approval date: 2016-09-28. Type: New approval. Active ingredient(s): Budesonide.
Notes: A drug with a new route of administration indicated for the treatment of mild to moderately active Crohn's disease.